PMDA approval: Mefeego Pack
Applicant: Linepharma K.K. Approval date: 2023-04-28. Type: New approval. Active ingredient(s): Mifepristone Misoprostol.
Notes: A drug with a new active ingredient and a new route of administration indicated for medical abortion within 63 days of gestation (9 weeks and 0 days of gestation) in women whose intrauterine pregnancy was confirmed.